PMDA approval: NovoSeven HI for Intravenous Injection 1 mg NovoSeven HI for Intravenous Injection 2 mg NovoSeven HI for Intravenous Injection 5 mg
Applicant: Novo Nordisk Pharma Ltd. Approval date: 2013-05-16. Type: Partial change. Active ingredient(s): Eptacog alfa (activated) (genetical recombination).
Notes: Drugs with a new dosage to add single-dose administration for the prevention of bleeding in patients with congenital hemophilia who have inhibitors against blood coagulation factor VIII or IX. [Public knowledge-based application after PAFSC's preliminary assessment]